PMDA approval: Latuda Tablets 20 mg Latuda Tablets 40 mg Latuda Tablets 60 mg Latuda Tablets 80 mg
Applicant: Sumitomo Dainippon Pharma Co., Ltd. Approval date: 2020-03-25. Type: New approval. Active ingredient(s): Lurasidone hydrochloride.
Notes: Drugs with a new active ingredient indicated for the treatment of schizophrenia and the improvement of depressive symptoms in patients with bipolar disorder.